PMDA approval: Agrylin Capsules 0.5 mg
Applicant: Shire Japan KK. Approval date: 2014-09-26. Type: New approval. Active ingredient(s): Anagrelide hydrochloride hydrate.
Notes: A drug with a new active ingredient indicated for the treatment of essential thrombocythemia. [Orphan drug]